PMDA approval: Herceptin Intravenous Infusion 60 Herceptin Intravenous Infusion 150
Applicant: Chugai Pharmaceutical Co., Ltd. Approval date: 2011-03-10. Type: Partial change. Active ingredient(s): Trastuzumab (genetical recombination).
Notes: Drugs with a new additional indication and a new dosage for the treatment of unresectable advanced or recurrent gastric cancer with HER2 overexpression. [Priority review]